PMDA approval: Adtralza S.C. Injection 150 mg Syringe
Applicant: Leo Pharma K.K. Approval date: 2022-12-23. Type: New approval. Active ingredient(s): Tralokinumab (genetical recombination).
Notes: A drug with a new active ingredient indicated for the treatment of atopic dermatitis in patients who have not responded sufficiently to conventional treatments.